PMDA approval: Sandostatin LAR for I.M. Injection 10 mg Sandostatin LAR for I.M. Injection 20 mg Sandostatin LAR for I.M. Injection 30 mg
Applicant: Novartis Pharma K.K. Approval date: 2011-11-25. Type: Partial change. Active ingredient(s): Octreotide acetate.
Notes: Drugs with a new additional indication and a new dosage for the treatment of gastrointestinal neuroendocrine tumor. [Public knowledge-based application after PAFSC's preliminary assessment]